PMDA approval: Alesion Eyelid Cream 0.5%
Applicant: Santen Pharmaceutical Co., Ltd. Approval date: 2024-03-26. Type: New approval. Active ingredient(s): Epinastine hydrochloride.
Notes: A drug in a new dosage form indicated for the treatment of allergic conjunctivitis.